PMDA approval: Gemzar Injection 200 mg Gemzar Injection 1 g
Applicant: Eli Lilly Japan K.K. Approval date: 2010-02-05. Type: Partial change. Active ingredient(s): Gemcitabine hydrochloride.
Notes: Drugs with a new additional indication and a new dosage for the treatment of inoperable or recurrent breast cancer.